PMDA approval: Bevacizumab BS Intravenous Infusion 100 mg "Nichi-Iko" Bevacizumab BS Intravenous Infusion 400 mg "Nichi-Iko"
Applicant: Nichi-Iko Pharmaceutical Co., Ltd. Approval date: 2022-01-20. Type: New approval. Active ingredient(s): Bevacizumab (genetical recombination) [bevacizumab biosimilar 3].
Notes: Follow-on biologics indicated for the treatment of unresectable advanced or recurrent colon or rectal cancer and unresectable advanced or recurrent non-squamous non-small cell lung cancer.